PMDA approval: Lunabell Tablets ULD
Applicant: Nobelpharma Co., Ltd. Approval date: 2022-03-11. Type: Partial change. Active ingredient(s): Norethisterone/ Ethinylestradiol.
Notes: A drug with a new indication and a new dosage for adjusting the start timing of controlled ovarian stimulation in assisted reproductive technology. [Public knowledge-based application]